PMDA approval: Azilva Tablets 10 mg Azilva Tablets 20 mg Azilva Granules 1%
Applicant: Takeda Pharmaceutical Company Limited. Approval date: 2024-08-28. Type: Partial change. Active ingredient(s): Azilsartan.
Notes: Drugs with a new additional pediatric dosage (aged 2 years to 5 years) indicated for the treatment of hypertension.